PMDA approval: E Keppra Tablets 250 mg E Keppra Tablets 500 mg E Keppra Dry Syrup 50% E Keppra for I.V. Infusion 500 mg
Applicant: UCB Japan Co., Ltd. Approval date: 2015-02-20. Type: Partial change. Active ingredient(s): Levetiracetam.
Notes: Drugs with a revised indication and a new dosage for the treatment of partial seizure in patients with epilepsy (including secondary generalized seizure).